PMDA approval: Vimpat Tablets 50 mg Vimpat Tablets 100 mg
Applicant: UCB Japan Co., Ltd. Approval date: 2016-07-04. Type: New approval. Active ingredient(s): Lacosamide.
Notes: Drugs with a new active ingredient indicated for the use as an adjunctive therapy with other antiepileptic drugs to treat partial seizures (including secondary generalized seizures) in patients with epilepsy who have not responded sufficiently to other antiepileptic drugs.